PMDA approval: Libtayo I.V. Infusion 350 mg
Applicant: Regeneron Japan KK. Approval date: 2025-09-19. Type: Partial change. Active ingredient(s): Cemiplimab (genetical recombination).
Notes: A drug with a new indication for the treatment of unresectable advanced or recurrent non-small-cell lung cancer.